PMDA approval: Isentress Tablets 400 mg
Applicant: Banyu Pharmaceutical Co., Ltd. Approval date: 2008-06-24. Type: New approval. Active ingredient(s): Raltegravir potassium.
Notes: A drug containing a new active ingredient indicated for the treatment of HIV infection. [Orphan drug]